PMDA approval: Polivy for Intravenous Infusion 140 mg Polivy for Intravenous Infusion 30 mg
Applicant: Chugai Pharmaceutical Co., Ltd. Approval date: 2021-03-23. Type: New approval. Active ingredient(s): Polatuzumab vedotin (genetical recombination).
Notes: Drugs with a new active ingredient indicated for the treatment of relapsed or refractory diffuse large B-cell lymphoma. [Orphan drug]